PMDA approval: Imbruvica Capsules 140 mg
Applicant: Janssen Pharmaceutical K.K. Approval date: 2022-12-23. Type: Partial change. Active ingredient(s): Ibrutinib.
Notes: A drug with new indications for the treatment of Waldenström's macroglobulinemia and lymphoplasmacytic lymphoma. [Orphan drug]